PMDA approval: Xarelto Tablets 10 mg Xarelto Tablets 15 mg
Applicant: Bayer Yakuhin, Ltd. Approval date: 2015-09-24. Type: Partial change. Active ingredient(s): Rivaroxaban.
Notes: Drugs with a new additional indication and a new dosage for the treatment and prevention of recurrence of deep vein thrombosis and pulmonary thromboembolism.